PMDA approval: Altuviiio Intravenous 250 Altuviiio Intravenous 500 Altuviiio Intravenous 1000 Altuviiio Intravenous 2000 Altuviiio Intravenous 3000 Altuviiio Intravenous 4000
Applicant: Sanofi K.K. Approval date: 2023-09-25. Type: New approval. Active ingredient(s): Efanesoctocog alfa (genetical recombination).
Notes: Drugs with a new active ingredient indicated for the control of bleeding tendency in patients with blood coagulation factor VIII deficiency.